PMDA approval: Zicthoru Tapes 75 mg
Applicant: Hisamitsu Pharmaceutical Co., Inc. Approval date: 2021-03-23. Type: New approval. Active ingredient(s): Diclofenac sodium.
Notes: A drug with a new indication in a new dosage form for analgesia in various types of cancer.